PMDA approval: Glufast Tab. 5 mg Glufast Tab. 10 mg
Applicant: Kissei Pharmaceutical Co., Ltd. Approval date: 2009-02-23. Type: Partial change. Active ingredient(s): Mitiglinide calcium hydrate.
Notes: Drugs with a new additional indication for the treatment of postprandial glucose excursions in Type 2 diabetes mellitus in patients who have not benefited sufficiently from taking thiazolidines in conjunction with dietary and exercise regimens.